PMDA approval: Vfend Tablets 50 mg Vfend Tablets 200 mg Vfend for Intravenous Use 200 mg Vfend Dry Syrup 2800 mg
Applicant: Pfizer Japan Inc. Approval date: 2015-08-24. Type: Partial change. Active ingredient(s): Voriconazole.
Notes: Drugs with a new additional indication and new dosages for the prophylaxis of deep mycosis in patients after hematopoietic stem cell transplantation.